PMDA approval: Oxarol Ointment 25 μg/g Oxarol Lotion 25 μg/g
Applicant: Chugai Pharmaceutical Co., Ltd. Approval date: 2008-11-25. Type: Partial change. Active ingredient(s): Maxacalcitol.
Notes: Drugs with a new indication for the treatment of palmoplantar pustulosis.